PMDA approval: Cylocide N Injection 400 mg Cylocide N Injection 1 g
Applicant: Nippon Shinyaku Co., Ltd. Approval date: 2023-05-25. Type: Partial change. Active ingredient(s): Cytarabine.
Notes: Drugs with a new indication and a new dosage for the treatment of acute leukemia. [Public knowledge-based application, Expedited review]